PMDA approval: Harvoni Combination Tablets
Applicant: Gilead Sciences, Inc. Approval date: 2015-07-03. Type: New approval. Active ingredient(s): Ledipasvir acetonate/Sofosbuvir.
Notes: A new combination drug with new active ingredients indicated for the improvement of viremia in patients with chronic hepatitis C or compensated cirrhosis type C in serogroup 1 (genotype 1). [Priority review]